PMDA approval: Zithromac Intravenous Use 500 mg Zithromac Tablets 250 mg
Applicant: Pfizer Japan Inc. Approval date: 2012-06-22. Type: Partial change. Active ingredient(s): Azithromycin hydrate.
Notes: Drugs with a new additional indication and a new dosage for the treatment of pelvic inflammatory diseases.